PMDA approval: Skyrizi 75 mg for S.C. Injection Syringe 0.83 mL
Applicant: AbbVie GK. Approval date: 2019-03-26. Type: New approval. Active ingredient(s): Risankizumab (genetical recombination).
Notes: A drug with a new active ingredient indicated for the treatment of plaque psoriasis, psoriatic arthritis, pustular psoriasis and erythrodermic psoriasis in patients who have not responded sufficiently to conventional therapies.